PMDA approval: Demser Capsules 250 mg
Applicant: Ono Pharmaceutical Co., Ltd. Approval date: 2019-01-08. Type: New approval. Active ingredient(s): Metirosine.
Notes: A drug with a new active ingredient indicated for the improvement of the status of excessive catecholamine secretion in patients with pheochromocytoma. [Orphan drug]